PMDA approval: Abraxane I.V. Infusion 100 mg
Applicant: Taiho Pharmaceutical Co., Ltd. Approval date: 2014-12-18. Type: Partial change. Active ingredient(s): Paclitaxel.
Notes: A drug with a new additional indication and a new dosage for the treatment of unresectable pancreatic cancer. [Priority review]